PMDA approval: E Keppra Tablets 250 mg E Keppra Tablets 500 mg E Keppra Dry Syrup 50% E Keppra for I.V. Infusion 500 mg
Applicant: UCB Japan Co., Ltd. Approval date: 2016-02-29. Type: Partial change. Active ingredient(s): Levetiracetam.
Notes: Drugs with a new additional indication for use as an adjunctive therapy with other antiepileptic drugs to treat tonic-clonic seizures in patients with epilepsy who have not responded sufficiently to other antiepileptic drugs.